PMDA approval: Selara Tablets 25 mg Selara Tablets 50 mg Selara Tablets 100 mg
Applicant: Pfizer Japan Inc. Approval date: 2007-07-31. Type: New approval. Active ingredient(s): Eplerenone.
Notes: Drugs containing a new active ingredient indicated for the treatment of hypertension.